PMDA approval: Doptelet Tablets 20 mg
Applicant: Swedish Orphan Biovitrum Japan Co., Ltd. Approval date: 2025-08-25. Type: Partial change. Active ingredient(s): Avatrombopag maleate.
Notes: A drug with a new indication and a new dosage for the treatment of persistent and chronic immune thrombocytopenia.